PMDA approval: Ondexxya for Intravenous Injection 200 mg
Applicant: Alexion Pharma G.K. Approval date: 2022-03-28. Type: New approval. Active ingredient(s): Andexanet alfa (genetical recombination).
Notes: A drug with a new active ingredient indicated for the reversal of anticoagulant effect in life-threatening or uncontrolled bleeding in patients treated with a direct-acting factor Xa inhibitor (apixaban, rivaroxaban, or edoxaban tosilate hydrate). [Orphan drug]